PMDA approval: Dalacin S Injection 300 mg Dalacin S Injection 600 mg
Applicant: Pfizer Japan Inc. Approval date: 2014-02-21. Type: Partial change. Active ingredient(s): Clindamycin phosphate.
Notes: Drugs with a new additional indication for cellulitis around the jaw bone and jaw inflammation. [Public knowledge-based application after PAFSC's preliminary assessment]